PMDA approval: Topina Tablets 25 mg Topina Tablets 50 mg Topina Tablets 100 mg
Applicant: Kyowa Hakko Kirin Co., Ltd. Approval date: 2013-11-22. Type: Partial change. Active ingredient(s): Topiramate.
Notes: Drugs with a new additional pediatric dosage. These drugs are indicated for use as an adjunctive therapy with other antiepileptic drugs to treat partial seizures (including secondary generalized seizures) in patients with epilepsy who have not responded sufficiently to other antiepileptic drugs.